PMDA approval: Orfadin Capsules 2 mg Orfadin Capsules 5 mg Orfadin Capsules 10 mg
Applicant: Astellas Pharma Inc. Approval date: 2014-12-26. Type: New approval. Active ingredient(s): Nitisinone.
Notes: Drugs with a new active ingredient indicated for the treatment of type I hypertyrosinemia.